PMDA approval: Nexium Capsules 10 mg
Applicant: AstraZeneca K.K. Approval date: 2011-07-01. Type: New approval. Active ingredient(s): Esomeprazole magnesium hydrate.
Notes: A drug with a new active ingredient indicated for the treatment of gastric ulcer, duodenal ulcer, anastomotic ulcer, reflux esophagitis, non-erosive reflux disease and Zollinger-Ellison syndrome, prevention of recurrence of gastric ulcer or duodenal ulcer in patients treated with nonsteroidal antiinflammatory drugs, and aid to eradication of Helicobacter pylori in patients with gastric ulcer, duodenal ulcer, gastric MALT lymphoma, idiopathic thrombocytopenic purpura, and stomach after endoscopic treatment for early gastric cancer. Nexium Capsules 20 mg (AstraZeneca K.K.) Approval Esomeprazole magnesium hydrate A drug with a new active ingredient indicated for the treatment of gastric ulcer, duodenal ulcer, anastomotic ulcer, reflux esophagitis and Zollinger-Ellison syndrome, prevention of recurrence of gastric ulcer or duodenal ulcer in patients treated with nonsteroidal antiinflammatory drugs, and aid to eradication of Helicobacter pylori in patients with gastric ulcer, duodenal ulcer, gastric MALT lymphoma, idiopathic thrombocytopenic purpura, and stomach after endoscopic treatment for early gastric cancer.